PMDA approval: Somatuline 60 mg for s.c. Injection Somatuline 90 mg for s.c. Injection Somatuline 120 mg for s.c. Injection
Applicant: Teijin Pharma Limited. Approval date: 2020-12-25. Type: Partial change. Active ingredient(s): Lanreotide acetate.
Notes: Drugs with a new indication for the treatment of thyroid-stimulating hormone secreting pituitary tumour. [Orphan drug]